PMDA approval: Bavencio Injection 200 mg
Applicant: Merck Biopharma Co., Ltd. Approval date: 2021-02-24. Type: Partial change. Active ingredient(s): Avelumab (genetical recombination).
Notes: A drug with a new indication for the maintenance therapy of unresectable urothelial carcinoma in patients who have received chemotherapy. [Priority review]